PMDA approval: Shingrix for Intramuscular Injection
Applicant: Japan Vaccine Co., Ltd. Approval date: 2018-03-23. Type: New approval. Active ingredient(s): Freeze-dried recombinant herpes zoster vaccine (prepared from Chinese hamster ovary cells).
Notes: A drug with a new active ingredient indicated for the prevention of herpes zoster.